PMDA approval: Tecentriq Intravenous Infusion 1200 mg
Applicant: Chugai Pharmaceutical Co., Ltd. Approval date: 2018-12-21. Type: Partial change. Active ingredient(s): Atezolizumab (genetical recombination).
Notes: A drug with a new dosage indicated for the treatment of unresectable advanced or recurrent non-small-cell lung cancer. [Priority review]